PMDA approval: Rekovelle Pen for S.C. Injection 12 µg Rekovelle Pen for S.C. Injection 36 µg Rekovelle Pen for S.C. Injection 72 µg
Applicant: Ferring Pharmaceuticals Co., Ltd. Approval date: 2021-03-23. Type: New approval. Active ingredient(s): Follitropin delta (genetical recombination).
Notes: Drugs with a new active ingredient indicated for controlled ovarian stimulation in assisted reproductive technology.